PMDA approval: Valixa Tablets 450 mg
Applicant: Mitsubishi Tanabe Pharma Corporation. Approval date: 2016-08-26. Type: Partial change. Active ingredient(s): Valganciclovir hydrochloride.
Notes: A drug with a new additional indication and a new dosage for the prevention of cytomegalovirus disease in organ transplant patients (excluding hematopoietic stem cell transplantation). [Public knowledge-based application after PAFSC's preliminary assessment]